PMDA approval: Rituxan Injection 10 mg/mL
Applicant: Zenyaku Kogyo Co., Ltd. Approval date: 2017-06-26. Type: Partial change. Active ingredient(s): Rituximab (genetical recombination).
Notes: A drug with a new additional indication and a new dosage for the treatment of chronic idiopathic thrombocytopenic purpura. [Public knowledge-based application after preliminary assessment by the Pharmaceutical Affairs and Food Sanitation Council (PAFSC)]